PMDA approval: Nobelzin Capsules 25 mg Nobelzin Capsules 50 mg
Applicant: Nobelpharma Co., Ltd. Approval date: 2008-01-25. Type: New approval. Active ingredient(s): Zinc acetate dihydrate.
Notes: Drugs containing a new active ingredient indicated for the treatment of Wilson’s disease (hepatolenticular degeneration). [Orphan drug]